PMDA approval: Micombi Combination Tablets AP
Applicant: Nippon Boehringer Ingelheim Co., Ltd. Approval date: 2009-04-22. Type: New approval. Active ingredient(s): Telmisartan/ hydrochlorothiazide.
Notes: New combination drugs indicated for the treatment of hypertension. 10 Micombi Combination Tablets BP (Nippon Boehringer Ingelheim Co., Ltd.) Approval